PMDA approval: Tecentriq Intravenous Infusion 1200 mg Tecentriq Intravenous Infusion 840 mg
Applicant: Chugai Pharmaceutical Co., Ltd. Approval date: 2025-02-20. Type: Partial change. Active ingredient(s): Atezolizumab (genetical recombination).
Notes: Drugs with a new indication and a new dosage for the treatment of unresectable alveolar soft part sarcoma.